PMDA approval: Cyramza Injection 100 mg Cyramza Injection 500 mg
Applicant: Eli Lilly Japan K.K. Approval date: 2019-06-18. Type: Partial change. Active ingredient(s): Ramucirumab (genetical recombination).
Notes: Drugs with a new indication for the treatment of patients with unresectable hepatocellular carcinoma whose serum AFP level is greater than 400 ng/mL and conditions have progressed after cancer chemotherapy.